PMDA approval: Izervay for Intravitreal Injection 20 mg/mL
Applicant: Astellas Pharma Inc. Approval date: 2025-09-19. Type: New approval. Active ingredient(s): Avacincaptad pegol sodium.
Notes: A drug with a new active ingredient indicated for slowing the progression of geographic atrophy secondary to age-related macular degeneration. [Conditional approval]